PMDA approval: Vasolan Tablets 40 mg
Applicant: Eisai Co., Ltd. Approval date: 2011-05-20. Type: Partial change. Active ingredient(s): Verapamil hydrochloride.
Notes: A drug with a new additional indication and a new dosage for the treatment of tachyarrhythmia in pediatric patients (atrial fibrillation, atrial flutter, paroxysmal supraventricular tachycardia). [Public knowledge-based application after PAFSC's preliminary assessment]